PMDA approval: Pentcillin for Injection 1 g Pentcillin for Injection 2 g Pentcillin Intravenous Injection 1 g Bag Pentcillin Intravenous Injection 2 g Bag
Applicant: Toyama Chemical Co., Ltd. Approval date: 2015-03-20. Type: New approval. Active ingredient(s): Piperacillin sodium.
Notes: Drugs with a new dosage indicated for the treatment of sepsis, acute bronchitis, pneumonia, lung abscess, thoracic empyema, secondary infection of chronic respiratory disease, cystitis, pyelonephritis, cholecystitis, choledochitis, bartholinitis, intrauterine infection, uterine adnexitis, parametritis, and purulent meningitis.